PMDA approval: Stivarga Tablets 40 mg
Applicant: Bayer Yakuhin, Ltd. Approval date: 2017-06-26. Type: Partial change. Active ingredient(s): Regorafenib hydrate.
Notes: A drug with a new additional indication for the treatment of unresectable hepatocellular carcinoma which has progressed after cancer chemotherapy. [Priority review]